PMDA approval: (1) Cellcept Capsules 250 (2) Cellcept Powder for Oral Suspension 31.8% (3) Mycophenolate Mofetil Capsules 250 mg [VTRS]
Applicant: [1] [2] Chugai Pharmaceutical Co., Ltd., [3] Viatris Healthcare G.K. Approval date: 2025-09-19. Type: Partial change. Active ingredient(s): Mycophenolate mofetil.
Notes: Drugs with a new indication and a new dosage for the treatment of refractory nephrotic syndrome (for use frequent relapsing or steroid-dependent nephrotic syndrome). [Public knowledge-based application after preliminary assessment by the Pharmaceutical Affairs and Food Sanitation Council (PAFSC)]